PMDA approval: Omjjara Tablets 100 mg Omjjara Tablets 150 mg Omjjara Tablets 200 mg
Applicant: GlaxoSmithKline K.K. Approval date: 2024-06-24. Type: New approval. Active ingredient(s): Momelotinib hydrochloride hydrate.
Notes: Drugs with a new active ingredient indicated for the treatment of myelofibrosis.